PMDA approval: Amvuttra Subcutaneous Injection 25 mg Syringe
Applicant: Alnylam Japan, K.K. Approval date: 2025-06-24. Type: Partial change. Active ingredient(s): Vutrisiran sodium.
Notes: A drug with a new indication for the treatment of transthyretin cardiac amyloidosis (wild-type and hereditary). [Orphan drug]